PMDA approval: Kenketsu Venilon-I for Intravenous Injection 500 mg Kenketsu Venilon-I for Intravenous Injection 1000 mg Kenketsu Venilon-I for Intravenous Injection 2500 mg Kenketsu Venilon-I for Intravenous Injection 5000 mg
Applicant: KM Biologics Co., Ltd. Approval date: 2019-12-20. Type: Partial change. Active ingredient(s): Freeze-dried sulfonated human normal immunoglobulin.
Notes: Drugs with a new additional indication and a new dosage for the treatment of acute-phase optic neuritis (for use when steroid drugs are not sufficiently effective). [Orphan drug]